PMDA approval: Bosulif Tablets 100 mg
Applicant: Pfizer Japan Inc. Approval date: 2014-09-26. Type: New approval. Active ingredient(s): Bosutinib hydrate.
Notes: A drug with a new active ingredient indicated for the treatment of chronic myelogenous leukemia with resistance or intolerance to prior drug therapies. [Orphan drug]